PMDA approval: Pemetrexed for I.V. Infusion 100 mg “NK” Pemetrexed for I.V. Infusion 500 mg “NK” Pemetrexed for I.V. Infusion 800 mg “NK” Pemetrexed Solution for I.V. Infusion 100 mg “NK” Pemetrexed Solution for I.V. Infusion 500 mg “NK” Pemetrexed Solution for I.V. Infusion 800 mg “NK”
Applicant: Nippon Kayaku Co., Ltd. Approval date: 2024-08-28. Type: Partial change. Active ingredient(s): Pemetrexed sodium hemipentahydrate.
Notes: Drugs with a new dosage for the preoperative adjuvant treatment for non-squamous non-small cell lung cancer.